PMDA approval: Thaled Capsules 25 Thaled Capsules 50 Thaled Capsules 100
Applicant: Fujimoto Pharmaceutical Corporation. Approval date: 2021-02-24. Type: Partial change. Active ingredient(s): Thalidomide.
Notes: Drugs with a new indication and a new dosage for the treatment of Crow-Fukase (polyneuropathy, organomegaly, endocrinopathy, M-protein, and skin changes [POEMS]) syndrome. [Orphan drug]